PMDA approval: Lupkynis Capsules 7.9 mg
Applicant: Otsuka Pharmaceutical Co., Ltd. Approval date: 2024-09-24. Type: New approval. Active ingredient(s): Voclosporin.
Notes: A drug with a new active ingredient indicated for the treatment of lupus nephritis.